PMDA approval: Levofolinate for I.V. Infusion 25 mg “Yakult” Levofolinate for I.V. Infusion 100 mg “Yakult”
Applicant: Yakult Honsha Co., Ltd. Approval date: 2013-12-20. Type: Partial change. Active ingredient(s): Levofolinate calcium.
Notes: Drugs with a new additional indication and a new dosage for the treatment of unresectable pancreatic cancer. [Priority review], [Expedited review]